PMDA approval: Vimpat Tablets 50 mg Vimpat Tablets 100 mg
Applicant: UCB Japan Co., Ltd. Approval date: 2017-08-25. Type: Partial change. Active ingredient(s): Lacosamide.
Notes: Drugs with a revised indication for the treatment of partial seizures (including secondary generalized seizures) in patients with epilepsy.